PMDA approval: Alimta Injection 100 mg Alimta Injection 500 mg
Applicant: Eli Lilly Japan K.K. Approval date: 2009-05-20. Type: New approval. Active ingredient(s): Pemetrexed sodium hydrate.
Notes: Drugs with a new additional indication and a new dosage in an additional dosage form (Alimta Injection 100 mg) indicated for the treatment of unresectable advanced or recurrent non-small cell lung cancer.